PMDA approval: Bonviva Tablet 100 mg
Applicant: Chugai Pharmaceutical Co., Ltd. Approval date: 2016-01-22. Type: New approval. Active ingredient(s): Ibandronate sodium hydrate.
Notes: A drug with a new route of administration indicated for the treatment of osteoporosis.